PMDA approval: Yervoy Injection 20 mg Yervoy Injection 50 mg
Applicant: Bristol-Myers Squibb K.K. Approval date: 2025-06-24. Type: Partial change. Active ingredient(s): Ipilimumab (genetical recombination).
Notes: Drugs with a new indication and a new dosage for the treatment of unresectable hepatocellular carcinoma.